PMDA approval: Cardiolite Injection Daiichi Change Technetium (99mTc) hexakis (2-methoxy-isobutyl isonitrile) Drugs with a new additional indication for diagnostic localization of hyperparathyroidism in parathyroid scintigraphy. Cardiolite Daiichi
Applicant: Fujifilm RI Pharma Co., Ltd. Approval date: 2010-02-05. Type: Partial change. Active ingredient(s): Tetrakis (2-methoxy-isobutyl isonitrile) copper (I) tetrafluoroborate.